PMDA approval: Nexviazyme for I.V. Infusion 100 mg
Applicant: Sanofi K.K. Approval date: 2021-09-27. Type: New approval. Active ingredient(s): Avalglucosidase alfa (genetical recombination).
Notes: A drug with a new active ingredient indicated for the treatment of Pompe disease. [Orphan drug]